PMDA approval: (1) Relvar 50 Ellipta 14 dose for Pediatric (2) Relvar 50 Ellipta 30 dose for Pediatric (3) Relvar 100 Ellipta 14 dose (4) Relvar 100 Ellipta 30 dose
Applicant: GlaxoSmithKline K.K. Approval date: 2024-06-24. Type: New approval. Active ingredient(s): Fluticasone furoate/Vilanterol trifenatate.
Notes: (1) (2) New combination drugs with a new additional pediatric dosage indicated for the treatment of bronchial asthma (when a combination treatment of an inhaled steroid and a long-acting beta-2 agonist is needed). (3) (4) Drugs with a new additional pediatric dosage indicated for the treatment of bronchial asthma (when a combination treatment of an inhaled steroid and a long-acting beta-2 agonist is needed).